PMDA approval: Forxiga Tablets 5 mg Forxiga Tablets 10 mg
Applicant: Bristol-Myers K.K. Approval date: 2014-03-24. Type: New approval. Active ingredient(s): Dapagliflozin propylene glycolate hydrate.
Notes: Drugs with a new active ingredient indicated for the treatment of type 2 diabetes mellitus.